PMDA approval: (1) Sogroya Subcutaneous Injection 5 mg (1) Sogroya Subcutaneous Injection 10 mg (2) Sogroya Subcutaneous Injection 15 mg
Applicant: Novo Nordisk Pharma Ltd. Approval date: 2023-06-26. Type: Partial change. Active ingredient(s): Somapacitan (genetical recombination).
Notes: (1) Drugs with a new indication, a new dosage, and other characteristics for the treatment of short stature due to growth hormone deficiency prior to epiphyseal closure. (2) A drug with a new indication and a new dosage in an additional dosage form for the treatment of short stature due to growth hormone deficiency prior to epiphyseal closure.